PMDA approval: Paxil Tablets 10 mg Paxil Tablets 20 mg
Applicant: GlaxoSmithKline K.K. Approval date: 2009-10-16. Type: Partial change. Active ingredient(s): Paroxetine hydrochloride hydrate.
Notes: Drugs with a new additional indication and a new dosage for the treatment of social anxiety disorder.